PMDA approval: Yervoy Injection 50 mg
Applicant: Bristol-Myers K.K. Approval date: 2015-07-03. Type: New approval. Active ingredient(s): Ipilimumab (genetical recombination).
Notes: A drug with a new active ingredient indicated for the treatment of unresectable melanoma. [Orphan drug]